PMDA approval: Xeljanz Tablets 5 mg
Applicant: Pfizer Japan Inc. Approval date: 2013-03-25. Type: New approval. Active ingredient(s): Tofacitinib citrate.
Notes: A drug with a new active ingredient for the treatment of rheumatoid arthritis in patients who have not sufficiently responded to conventional treatments.